PMDA approval: Quviviq Tablets 25 mg Quviviq Tablets 50 mg
Applicant: Nexera Pharma Co., Ltd. Approval date: 2024-09-24. Type: New approval. Active ingredient(s): Daridorexant hydrochloride.
Notes: Drugs with a new active ingredient indicated for the treatment of insomnia.